PMDA approval: Cyramza Injection 100 mg Cyramza Injection 500 mg
Applicant: Eli Lilly Japan K.K. Approval date: 2016-05-23. Type: Partial change. Active ingredient(s): Ramucirumab (genetical recombination).
Notes: Drugs with a new additional indication and a new dosage for the treatment of unresectable advanced or recurrent colorectal cancer.